PMDA approval: Careload LA Tablets 60 µg (Toray Industries, Inc.) Berasus LA Tablets 60 µg
Applicant: Kaken Pharmaceutical Co., Ltd. Approval date: 2007-10-19. Type: New approval. Active ingredient(s): Beraprost sodium.
Notes: Drugs with a new indication and dosage and in a new dosage form for the treatment of pulmonary arterial hypertension. [Priority review]